PMDA approval: Tin Colloid Tc-99m Kit
Applicant: Nihon Medi-Physics Co., Ltd. Approval date: 2009-09-18. Type: Partial change. Active ingredient(s): Technetium (99mTc) stannous colloid.
Notes: A drug with a new route of administration and a new indication for sentinel lymph node mapping in breast cancer and malignant melanoma. [Expedited review]